PMDA approval: Eylea Intravitreal Injection 40 mg/mL Eylea Intravitreal Injection Kit 40 mg/mL
Applicant: Bayer Yakuhin, Ltd. Approval date: 2020-03-25. Type: Partial change. Active ingredient(s): Aflibercept (genetical recombination).
Notes: Drugs with a new indication and a new dosage for the treatment of neovascular glaucoma. [Orphan drug]